PMDA approval: Brukinsa Capsules 80 mg
Applicant: BeiGene Japan GK. Approval date: 2024-12-27. Type: New approval. Active ingredient(s): Zanubrutinib.
Notes: A drug with a new active ingredient indicated for the treatment of chronic lymphocytic leukemia (including small lymphocytic lymphoma), Waldenström's macroglobulinemia, and lymphoplasmacytic lymphoma.